PMDA approval: Cedarcure Japanese Cedar Pollen Sublingual Tablets 2,000 JAU Cedarcure Japanese Cedar Pollen Sublingual Tablets 5,000 JAU
Applicant: Torii Pharmaceutical Co., Ltd. Approval date: 2017-09-27. Type: New approval. Active ingredient(s): Japanese cedar pollen extract bulk powder.
Notes: Drugs with a new active ingredient indicated for the treatment of Japanese cedar pollinosis (desensitization therapy).